PMDA approval: Adynovate Intravenous Kit 250 Adynovate Intravenous Kit 500 Adynovate Intravenous Kit 1000 Adynovate Intravenous Kit 1500 Adynovate Intravenous Kit 2000 Adynovate Intravenous Kit 3000
Applicant: Takeda Pharmaceutical Company Limited. Approval date: 2023-06-26. Type: Partial change. Active ingredient(s): Rurioctocog alfa pegol (genetical recombination).
Notes: Drugs with a new dosage indicated for the control of bleeding tendency in patients with blood coagulation factor VIII deficiency.